PMDA approval: Bisono Tape 4 mg Bisono Tape 8 mg
Applicant: Toa Eiyo Ltd. Approval date: 2013-06-28. Type: New approval. Active ingredient(s): Bisoprolol.
Notes: Drugs with a new active ingredient indicated for the treatment of essential hypertension (mild to moderate).